PMDA approval: Bepricor Tablets 50 mg Bepricor Tablets 100 mg
Applicant: Schering-plough K.K. Approval date: 2008-10-16. Type: Partial change. Active ingredient(s): Bepridil hydrochloride hydrate.
Notes: Drugs with a new indication for the treatment of persistent atrial fibrillation in patients in whom other antiarrhythmic drugs are contraindicated or who have not responded to other antiarrhythmic drugs. [Priority review]